PMDA approval: Bimzelx Syringe for S.C Injection 160 mg Bimzelx Autoinjector for S.C Injection 160 mg
Applicant: UCB Japan Co., Ltd. Approval date: 2023-12-22. Type: Partial change. Active ingredient(s): Bimekizumab (genetical recombination).
Notes: Drugs with a new indication and a new dosage for the treatment of psoriatic arthritis, ankylosing spondylitis, and non-radiographic axial spondyloarthritis in patients who have not sufficiently responded to conventional treatments.